PMDA approval: Miticure House Dust Mite Sublingual Tablets 3,300 JAU Miticure House Dust Mite Sublingual Tablets 10,000 JAU
Applicant: Torii Pharmaceutical Co., Ltd. Approval date: 2015-09-28. Type: New approval. Active ingredient(s): Dermatophagoides farinae extract, Dermatophagoides pteronyssinus extract.
Notes: Drugs with new active ingredients indicated for the allergen immunotherapy for house dust mite antigen-induced allergic rhinitis.